PMDA approval: Vabysmo Solution for Intravitreal Injection 120 mg/mL
Applicant: Chugai Pharmaceutical Co., Ltd. Approval date: 2024-03-26. Type: Partial change. Active ingredient(s): Faricimab (genetical recombination).
Notes: A drug with a new indication and a new dosage for the treatment of macular edema following retinal vein occlusion.